PMDA approval: Rheumatrex Capsules 2 mg (Pfizer Japan Inc.) Methotrexate Tablets 2 mg "Tanabe" (Mitsubishi Tanabe Pharma Corporation) Methotrexate Cap. 2 mg "Mylan" (Mylan Seiyaku Ltd.) Trexamette Capsules 2 mg (Shiono Chemical Co., Ltd.) Methotrexate Capsules 2 mg "Towa" (Towa Pharmaceutical Co., Ltd.) Metolate Tablets 2 mg (Santen Pharmaceutical Co., Ltd.) Methotrexate Cap. 2 mg "Sawai"
Applicant: Sawai Pharmaceutical Co., Ltd. Approval date: 2011-02-23. Type: Partial change. Active ingredient(s): Methotrexate.
Notes: Drugs with a revised indication and an expanded dosage for the treatment of rheumatoid arthritis (limitation of patients to be treated was abolished).